PMDA approval: Epadel EM Capsules 2 g
Applicant: Mochida Pharmaceutical Co., Ltd. Approval date: 2022-06-20. Type: New approval. Active ingredient(s): Ethyl Icosapentate.
Notes: A drug in a new dosage form indicated for the treatment of hyperlipidemia.